PMDA approval: Lucentis Solution for Intravitreal Injection 10 mg/mL
Applicant: Novartis Pharma K.K. Approval date: 2019-11-22. Type: Partial change. Active ingredient(s): Ranibizumab (genetical recombination).
Notes: A drug with a new additional indication and a new dosage for the treatment of retinopathy of prematurity. [Orphan drug]